PMDA approval: Prevenar 13 Suspension Liquid for Injection
Applicant: Pfizer Japan Inc. Approval date: 2013-06-18. Type: New approval. Active ingredient(s): Pneumococcal 13-valent conjugate vaccine adsorbed (mutated diphtheria CRM197 conjugate).
Notes: A drug with a new active ingredient indicated for the prophylaxis of pneumococcal invasive disease (serotypes 1, 3, 4, 5, 6A, 6B, 7F, 9V, 14, 18C, 19A, 19F, and 23F).